PMDA approval: Actos Tablets 15 Actos Tablets 30
Applicant: Takeda Pharmaceutical Company Limited. Approval date: 2008-12-22. Type: Partial change. Active ingredient(s): Pioglitazone hydrochloride.
Notes: Drugs with a new additional indication for the treatment of Type 2 diabetes mellitus (for use only in patients in whom treatment with biguanides in conjunction with dietary and exercise regimens is not sufficiently effective, and insulin resistance is suspected).